PMDA approval: Xofluza Tablets 10 mg Xofluza Tablets 20 mg
Applicant: Shionogi & Co., Ltd. Approval date: 2018-02-23. Type: New approval. Active ingredient(s): Baloxavir marboxil.
Notes: Drugs with a new active ingredient indicated for the treatment of influenza A or B virus infections. [SAKIGAKE designation]